PMDA approval: Takecab Tablets 10 mg Takecab Tablets 20 mg
Applicant: Takeda Pharmaceutical Company Limited. Approval date: 2014-12-26. Type: New approval. Active ingredient(s): Vonoprazan fumarate.
Notes: Drugs with a new active ingredient indicated for the treatment of gastric ulcer, duodenal ulcer, reflux esophagitis, prevention of recurrence of gastric or duodenal ulcer in patients treated with low-dose aspirin, prevention of recurrence of gastric or duodenal ulcer in patients treated with non-steroidal anti-inflammatory drugs (NSAIDs), and aid to eradication of Helicobacter pylori in patients with gastric ulcer, duodenal ulcer, gastric MALT lymphoma, idiopathic thrombocytopenic purpura, Helicobacter pylori gastritis, and stomach after endoscopic treatment for early gastric cancer.